PMDA approval: Synvisc Intraarticular Injection 2 mL
Applicant: Genzyme Japan K.K. Approval date: 2010-07-23. Type: New approval. Active ingredient(s): Sodium hyaluronate crosslinked polymer and sodium hyaluronate crosslinked polymer crosslinked with vinylsulfone.
Notes: A drug with a new active ingredient indicated for pain relief in patients with knee osteoarthritis who have not responded sufficiently to conservative non-drug treatment and oral drug treatment.